PMDA approval: Refixia I.V. Injection 500 Refixia I.V. Injection 1000 Refixia I.V. Injection 2000
Applicant: Novo Nordisk Pharma Ltd. Approval date: 2018-07-02. Type: New approval. Active ingredient(s): Nonacog beta pegol (genetical recombination).
Notes: Drugs with a new active ingredient indicated for the control of bleeding tendency in patients with coagulation factor IX deficiency.